PMDA approval: Rozerem Tablets 8 mg
Applicant: Takeda Pharmaceutical Company Limited. Approval date: 2010-04-16. Type: New approval. Active ingredient(s): Ramelteon.
Notes: A drug with a new active ingredient indicated for the improvement of difficulty with sleep onset in insomnia.